PMDA approval: Iomeron 350 Iomeron 350 Syringe
Applicant: Bracco-Eisai Co., Ltd. Approval date: 2008-05-20. Type: Partial change. Active ingredient(s): Iomeprol.
Notes: A drug with a new dosage (Iomeron 350) and a drug with a new dosage in a new dosage form (Iomeron 350 Syringe) indicated for use in angiocardiography, thoracic angiography, abdominal angiography, angiography of the extremities, intravenous digital angiography, arterial digital angiography, computed tomography, and intravenous urography.